PMDA approval: Ecclock Gel 5%
Applicant: Kaken Pharmaceutical Co., Ltd. Approval date: 2020-09-25. Type: New approval. Active ingredient(s): Sofpironium bromide.
Notes: A drug with a new active ingredient indicated for the treatment of primary axillary hyperhidrosis.